PMDA approval: Sivextro Tablets 200 mg Sivextro Intravenous Infusion 200 mg
Applicant: Bayer Yakuhin, Ltd. Approval date: 2018-03-23. Type: New approval. Active ingredient(s): Tedizolid phosphate.
Notes: Drugs with a new active ingredient indicated for the treatment of bacterial skin infections mainly involving the dermis and/or subcutaneous tissues, secondary bacterial infections of pre-existing skin ulcers and/or erosion, and secondary bacterial infections of trauma, burn, and surgical wounds.